PMDA approval: Ilaris for S.C. Injection 150 mg
Applicant: Novartis Pharma K.K. Approval date: 2016-12-19. Type: Partial change. Active ingredient(s): Canakinumab (genetical recombination).
Notes: A drug with new additional indications and a new dosage for the treatment of patients with familial Mediterranean fever who have not responded sufficiently to conventional therapies, TNF receptor-associated periodic syndrome, or hyper IgD syndrome (mevalonate kinase deficiency). [Orphan drug]